PMDA approval: Clexane Subcutaneous Injection Kit 2000 IU
Applicant: Sanofi-Aventis K.K. Approval date: 2008-01-25. Type: New approval. Active ingredient(s): Enoxaparin sodium.
Notes: A drug containing a new active ingredient indicated for the prevention of venous thromboembolism in patients who have undergone total hip replacement, total knee replacement and hip fracture surgery. [Priority review]